PMDA approval: Infliximab BS for I.V. Infusion 100 mg [Pfizer]
Applicant: Pfizer Japan Inc. Approval date: 2018-07-02. Type: New approval. Active ingredient(s): Infliximab (genetical recombination) [infliximab biosimilar 3].
Notes: Follow-on biologics indicated for the treatment of rheumatoid arthritis, psoriasis, Crohn’s disease, and ulcerative colitis.